PMDA approval: Rozex Gel 0.75%
Applicant: Galderma S.A. Approval date: 2014-12-26. Type: New approval. Active ingredient(s): Metronidazole.
Notes: A drug with a new route of administration indicated for disinfection/deodorisation of odorous fungating tumours.